PMDA approval: Imfinzi Injection 120 mg Imfinzi Injection 500 mg
Applicant: AstraZeneca K.K. Approval date: 2025-09-19. Type: Partial change. Active ingredient(s): Durvalumab (genetical recombination).
Notes: Drugs with a new indication and a new dosage for the preoperative and postoperative adjuvant treatment for non-small cell lung cancer and preoperative and postoperative adjuvant treatment for bladder cancer.